PMDA approval: Mundesine Capsule 100 mg
Applicant: Mundipharma K.K. Approval date: 2017-03-30. Type: New approval. Active ingredient(s): Forodesine hydrochloride.
Notes: A drug with a new active ingredient indicated for the treatment of relapsed or refractory peripheral T-cell lymphoma. [Orphan drug]